PMDA approval: Actemra 162 mg Syringe for SC Injection Actemra 162 mg Auto-injector for SC Injection
Applicant: Chugai Pharmaceutical Co., Ltd. Approval date: 2017-08-25. Type: Partial change. Active ingredient(s): Tocilizumab (genetical recombination).
Notes: Drugs with new additional indications and a new dosage for the treatment of Takayasu arteritis and giant cell arteritis in patients who have not responded sufficiently to conventional treatments. [Orphan drug]